PMDA approval: Alunbrig Tablets 30 mg Alunbrig Tablets 90 mg
Applicant: Takeda Pharmaceutical Company Limited. Approval date: 2021-01-22. Type: New approval. Active ingredient(s): Brigatinib.
Notes: Drugs with a new active ingredient indicated for the treatment of unresectable advanced/recurrent anaplastic lymphoma kinase (ALK)-positive non-small cell lung cancer.